PMDA approval: Allelock Tablets 2.5 Allelock Tablets 5 Allelock OD Tablets 2.5 Allelock OD Tablets 5
Applicant: Kyowa Hakko Kirin Co., Ltd. Approval date: 2010-07-23. Type: Partial change. Active ingredient(s): Olopatadine hydrochloride.
Notes: Drugs with a new additional pediatric dosage indicated for the treatment of allergic rhinitis, urticaria, and itching associated with skin disease (eczema/dermatitis and pruritus cutaneous).